PMDA approval: Venoglobulin IH 5% I.V. 2.5 g/50 mL
Applicant: Benesis Corporation. Approval date: 2010-05-13. Type: Partial change. Active ingredient(s): Polyethylene glycol treated human normal immunoglobulin.
Notes: A drug with a new dosage indicated for the treatment of hypogammaglobulinemia or agammaglobulinemia.